PMDA approval: Enoras Liquid for Enteral Use
Applicant: EN Otsuka Pharmaceutical Co., Ltd. Approval date: 2019-03-26. Type: New approval. Active ingredient(s): N/A for this combination drug.
Notes: A combination prescription drug with similar formulations indicated for tube feeding, especially for patients with long-term oral feeding difficulties. It also generally can be used for nutrient retention for postoperative patients.